PMDA approval: Acofide Tablets 100 mg
Applicant: Zeria Pharmaceutical Co., Ltd. Approval date: 2013-03-25. Type: New approval. Active ingredient(s): Acotiamide hydrochloride hydrate.
Notes: A drug with a new active ingredient indicated for the treatment of postprandial fullness, upper abdominal bloating, and early satiation in functional dyspepsia.